PMDA approval: Gemzar Injection 200 mg Gemzar Injection 1 g
Applicant: Eli Lilly Japan K.K. Approval date: 2011-02-23. Type: Partial change. Active ingredient(s): Gemcitabine hydrochloride.
Notes: Drugs with a new additional indication and a new dosage for the treatment of ovarian cancer which has progressed after cancer chemotherapy. [Public knowledge-based application after PAFSC's preliminary assessment]